PMDA approval: Carogra Tablets 120 mg
Applicant: EA Pharma Co., Ltd. Approval date: 2022-03-28. Type: New approval. Active ingredient(s): Carotegrast methyl.
Notes: A drug with a new active ingredient indicated for the treatment of moderate ulcerative colitis (for use only in patients who have not sufficiently responded to 5-aminosalicylic acid preparation).